PMDA approval: Emend Capsules 80 mg Emend Capsules 125 mg Emend Capsules Set
Applicant: Ono Pharmaceutical Co., Ltd. Approval date: 2012-06-22. Type: Partial change. Active ingredient(s): Aprepitant.
Notes: Drugs with a new additional pediatric dosage for patients of 12 years or older. These drugs are indicated for the treatment of gastrointestinal symptoms (nausea and vomiting) associated with administration of antineoplastic drugs (cisplatin, etc.) (including delayed phase).